PMDA approval: Plaquenil Tablets 200 mg
Applicant: Sanofi K.K. Approval date: 2015-07-03. Type: New approval. Active ingredient(s): Hydroxychloroquine sulfate.
Notes: A drug with a new active ingredient indicated for the treatment of cutaneous lupus erythematosus and systemic lupus erythematosus.